PMDA approval: Prezistanaive Tablets 400 mg
Applicant: Janssen Pharmaceutical K.K. Approval date: 2009-08-20. Type: New approval. Active ingredient(s): Darunavir ethanolate.
Notes: A drug with a new dosage indicated for the treatment of HIV infection. [Orphan drug]